PMDA approval: Tecentriq Intravenous Infusion 840 mg Tecentriq Intravenous Infusion 1200 mg
Applicant: Chugai Pharmaceutical Co., Ltd. Approval date: 2025-09-19. Type: Partial change. Active ingredient(s): Atezolizumab (genetical recombination).
Notes: Drugs with a new indication and a new dosage for the treatment of relapsed or refractory extranodal NK/T-cell lymphoma, nasal type.